PMDA approval: Tapenta Tablets 25 mg Tapenta Tablets 50 mg Tapenta Tablets 100 mg
Applicant: Janssen Pharmaceutical K.K. Approval date: 2014-03-24. Type: New approval. Active ingredient(s): Tapentadol hydrochloride.
Notes: Drugs with a new active ingredient indicated for management of moderate to severe pain in various types of cancer.